PMDA approval: Keytruda Injection 100 mg Keytruda Injection 20 mg
Applicant: MSD K.K. Approval date: 2016-12-19. Type: Partial change. Active ingredient(s): Pembrolizumab (genetical recombination).
Notes: Drugs with a new additional indication and a new dosage for the treatment of patients with PD-L1 - positive, unresectable, recurrent or advanced non-small cell lung cancer.